PMDA approval: Remicade for I.V. Infusion 100
Applicant: Mitsubishi Tanabe Pharma Corporation. Approval date: 2007-11-13. Type: Partial change. Active ingredient(s): Infliximab (genetically engineered).
Notes: A drug with a new indication and dosage for use as maintenance therapy in Crohn’s disease. [Orphan drug]